PMDA approval: Mitomycin 2 mg for Ophthalmic Topical Use
Applicant: Kyowa Kirin Co., Ltd. Approval date: 2022-12-23. Type: New approval. Active ingredient(s): Mitomycin C.
Notes: A drug with a new route of administration indicated for an adjunct to incisional glaucoma surgery.